PMDA approval: Neffy Nasal Spray 1 mg Neffy Nasal Spray 2 mg
Applicant: Alfresa Pharma Corporation. Approval date: 2025-09-19. Type: New approval. Active ingredient(s): Adrenaline.
Notes: Drugs with a new route of administration indicated for the adjunctive treatment for anaphylactic reactions caused by bee venom, foods, drugs, and others (for use only in individuals with a history of anaphylaxis or at a high risk of anaphylaxis).